PMDA approval: Zykadia Capsules 150 mg
Applicant: Novartis Pharma K.K. Approval date: 2016-03-28. Type: New approval. Active ingredient(s): Ceritinib.
Notes: A drug with a new active ingredient indicated for the treatment of unresectable advanced/relapsed anaplastic lymphoma kinase (ALK)-positive non-small-cell lung cancer with resistance or intolerance to crizotinib.